PMDA approval: Rapiacta 300 mg Bag for Intravenous Drip Infusion Rapiacta 150 mg Vial for Intravenous Drip Infusion
Applicant: Shionogi & Co., Ltd. Approval date: 2010-01-13. Type: New approval. Active ingredient(s): Peramivir hydrate.
Notes: Drugs with a new active ingredient indicated for the treatment of influenza A or B virus infections.